PMDA approval: Gonatropin 5000
Applicant: ASKA Pharmaceutical. Co., Ltd. Approval date: 2008-09-01. Type: Partial change. Active ingredient(s): [JP] Human chorionic gonadotrophin (extract from human urine).
Notes: A drug with a new route of administration and a new indication for induction of spermatogenesis in male hypogonadotropic hypogonadism. [Expedited review]